PMDA approval: Vyvdura Combination Subcutaneous Injection
Applicant: Argenx Japan K.K. Approval date: 2024-12-27. Type: Partial change. Active ingredient(s): Efgartigimod alfa (genetical recombination) and Vorhyaluronidase alfa (genetical recombination).
Notes: A drug with a new indication and a new dosage for the treatment of chronic inflammatory demyelinating polyneuropathy. [Orphan drug]